PMDA approval: Polivy for Intravenous Infusion 30 mg Polivy for Intravenous Infusion 140 mg
Applicant: Chugai Pharmaceutical Co., Ltd. Approval date: 2022-08-24. Type: Partial change. Active ingredient(s): Polatuzumab vedotin (genetical recombination).
Notes: Drugs with a new indication and a new dosage for the treatment of diffuse large B-cell lymphoma. [Orphan drug]